PMDA approval: Thaled Capsule 50 Thaled Capsule 100
Applicant: Fujimoto Pharmaceutical Corporation. Approval date: 2012-05-25. Type: Partial change. Active ingredient(s): Thalidomide.
Notes: Drugs with a new additional indication and a new dosage for the treatment of erythema nodosum leprosum. [Orphan drug]